PMDA approval: Pariet Tablets 5 mg Pariet Tablets 10 mg
Applicant: Eisai Co., Ltd. Approval date: 2014-12-26. Type: New approval. Active ingredient(s): Rabeprazole sodium.
Notes: A drug with a new additional indication and a new dosage in a newly-added dosage form, and a drug with a new additional indication and a new dosage for the prevention of recurrence of gastric ulcer or duodenal ulcer in patients treated with low-dose aspirin.